PMDA approval: Livalo Tablets 1 mg Livalo Tablets 2 mg Livalo Tablets 4 mg
Applicant: Kowa Company, Ltd. Approval date: 2013-02-28. Type: Partial change. Active ingredient(s): Pitavastatin calcium.
Notes: Drugs with a new dosage indicated for the treatment of hypercholesterolemia and familial hypercholesterolemia.